PMDA approval: Exelon Patch 4.5 mg Exelon Patch 9 mg Exelon Patch 13.5 mg Exelon Patch 18 mg (Novartis Pharma K.K.) Rivastach Patch 4.5 mg Rivastach Patch 9 mg Rivastach Patch 13.5 mg Rivastach Patch 18 mg
Applicant: Ono Pharmaceutical Co., Ltd. Approval date: 2011-04-22. Type: New approval. Active ingredient(s): Rivastigmine.
Notes: Drugs with a new active ingredient indicated for inhibition of progression of symptoms of dementia in mild and moderate Alzheimer's dementia.